PMDA approval: Gliadel for Intracerebral Implant 7.7 mg
Applicant: Nobelpharma Co., Ltd. Approval date: 2012-09-28. Type: New approval. Active ingredient(s): Carmustine.
Notes: A drug with a new active ingredient indicated for the treatment of malignant glioma. [Orphan drug]